PMDA approval: Taltz 80 mg Syringe for SC Injection Taltz 80 mg Auto-injector for SC Injection
Applicant: Eli Lilly Japan K.K. Approval date: 2019-11-22. Type: Partial change. Active ingredient(s): Ixekizumab (genetical recombination).
Notes: Drugs with a new additional indication and a new dosage for the treatment of ankylosing spondylitis in patients who have not responded sufficiently to conventional treatments.